PMDA approval: D Dry 2.75S
Applicant: Nikkiso Co., Ltd. Approval date: 2017-03-30. Type: New approval. Active ingredient(s): N/A for this combination drug.
Notes: A prescription drug with similar formulations to approved combination drugs used as perfusion fluids for hemodialysis in patients with chronic renal failure.